PMDA approval: Alaglio Divided Granules 1.5 g Alaglio Oral 1.5 g
Applicant: SBI Pharmaceuticals Co., Ltd. Approval date: 2024-09-24. Type: Partial change. Active ingredient(s): Aminolevulinic acid hydrochloride.
Notes: Drugs with a new dosage and other characteristics indicated for the visualization of tumor tissues of the non-muscle invasive bladder cancer in transurethral resection of bladder tumor. [Orphan drug]